PMDA approval: Lucentis Solution for Intravitreal Injection 2.3 mg/0.23 mL
Applicant: Novartis Pharma K.K. Approval date: 2009-01-21. Type: New approval. Active ingredient(s): Ranibizumab (genetical recombination).
Notes: A drug containing a new active ingredient indicated for treatment of age-related macular degeneration with concurrent choroidal neovascularization. [Orphan drug]